PMDA approval: Tenelia Tablets 20 mg
Applicant: Mitsubishi Tanabe Pharma Corporation. Approval date: 2012-06-29. Type: New approval. Active ingredient(s): Teneligliptin hydrobromide hydrate.
Notes: A drug with a new active ingredient indicated for the treatment of type 2 diabetes mellitus.